PMDA approval: Gadovist IV Inj. 1.0 mol/L 7.5 mL Gadovist IV Inj. 1.0 mol/L Syringe 5 mL Gadovist IV Inj. 1.0 mol/L Syringe 7.5 mL Gadovist IV Inj. 1.0 mol/L Syringe 10 mL
Applicant: Bayer Yakuhin, Ltd. Approval date: 2015-03-26. Type: New approval. Active ingredient(s): Gadobutrol.
Notes: Drugs with a new active ingredient indicated for visibility of brain/spinal cord and trunk/extremities in magnetic resonance computer tomographic imaging as a contrast agent.